PMDA approval: Farmorubicin for Injection 10 mg Farmorubicin for Injection 50 mg
Applicant: Pfizer Japan Inc. Approval date: 2013-11-22. Type: Partial change. Active ingredient(s): Epirubicin hydrochloride.
Notes: Drugs with a new additional dosage indicated for transcatheter arterial chemo-embolization (TACE) in hepatocellular carcinoma. [Public knowledge-based application]